PMDA approval: Vanflyta Tablets 17.7 mg Vanflyta Tablets 26.5 mg
Applicant: Daiichi Sankyo Company, Limited. Approval date: 2023-05-25. Type: Partial change. Active ingredient(s): Quizartinib hydrochloride.
Notes: Drugs with a new indication and a new dosage for the treatment of newly diagnosed FLT3-ITD mutation-positive acute myeloid leukemia. [Orphan drug]